PMDA approval: Lorbrena Tablets 25 mg Lorbrena Tablets 100 mg
Applicant: Pfizer Japan Inc. Approval date: 2018-09-21. Type: New approval. Active ingredient(s): Lorlatinib.
Notes: Drugs with a new active ingredient indicated for the treatment of unresectable or recurrent anaplastic lymphoma kinase (ALK)-positive non-small cell lung cancer with resistance or intolerance to ALK tyrosine kinase inhibitors. [Conditional early approval]